PMDA approval: Durotep MT Patch 2.1 mg Durotep MT Patch 4.2 mg Durotep MT Patch 8.4 mg Durotep MT Patch 12.6 mg Durotep MT Patch 16.8 mg
Applicant: Janssen Pharmaceutical K.K. Approval date: 2010-01-20. Type: Partial change. Active ingredient(s): Fentanyl.
Notes: Drugs with a new indication for analgesia of moderate to severe chronic pain which cannot be managed by treatments with non-opioid analgesics and weak opioid analgesics (for use only in patients who switch from an opioid analgesic).